PMDA approval: Revolade Tablets 12.5 mg Revolade Tablets 25 mg
Applicant: Novartis Pharma K.K. Approval date: 2023-12-22. Type: Partial change. Active ingredient(s): Eltrombopag olamine.
Notes: Drugs with a new additional pediatric dosage indicated for the treatment of aplastic anemia. [Orphan drug]